PMDA approval: Amenalief Tab. 200 mg
Applicant: Maruho Co., Ltd. Approval date: 2017-07-03. Type: New approval. Active ingredient(s): Amenamevir.
Notes: A drug with a new active ingredient indicated for the treatment of herpes zoster.